PMDA approval: Botox for Injection 50 Units Botox for Injection 100 Units
Applicant: GlaxoSmithKline K.K. Approval date: 2010-10-27. Type: Partial change. Active ingredient(s): Botulinum toxin type A.
Notes: Drugs with new additional indications and new dosages for the treatment of upper and lower limb spasticity.